PMDA approval: Olumiant Tablets 2 mg Olumiant Tablets 4 mg
Applicant: Eli Lilly Japan K.K. Approval date: 2021-04-23. Type: Partial change. Active ingredient(s): Baricitinib.
Notes: Drugs with a new indication and a new dosage for the treatment of pneumonia caused by SARS-CoV-2 (only for the patients requiring supplemental oxygen). [Priority review]